PMDA approval: Encruse 62.5 μg Ellipta 7 doses Encruse 62.5 μg Ellipta 30 doses
Applicant: GlaxoSmithKline K.K. Approval date: 2015-03-26. Type: New approval. Active ingredient(s): Umeclidinium bromide.
Notes: Other drugs with a new dosage indicated for the relief of symptoms secondary to airway obstructive disorder in chronic obstructive pulmonary disease (chronic bronchitis, emphysema).